PMDA approval: Arixtra Injection 1.5 mg Arixtra Injection 2.5 mg
Applicant: GlaxoSmithKline K.K. Approval date: 2008-05-20. Type: Partial change. Active ingredient(s): Fondaparinux sodium.
Notes: Drugs with a new indication for prophylaxis of venous thromboembolism in patients undergoing abdominal surgery who are at a high risk of developing venous thromboembolism. [Priority review]